PMDA approval: Cosentyx for S.C. Injection 150 mg Syringe Cosentyx for S.C. Injection 150 mg Pen
Applicant: Novartis Pharma K.K. Approval date: 2018-12-21. Type: Partial change. Active ingredient(s): Secukinumab (genetical recombination).
Notes: Drugs with a new additional indication and a new dosage for the treatment of ankylosing spondylitis in patients who have not responded sufficiently to conventional treatments.